PMDA approval: Zosyn for Intravenous Injection 2.25 Zosyn for Intravenous Injection 4.5
Applicant: Taiho Pharmaceutical Co., Ltd. Approval date: 2012-09-28. Type: Partial change. Active ingredient(s): Tazobactam/ piperacillin hydrate.
Notes: Drugs with new additional indications for the treatment of peritonitis, intraperitoneal abscess, cholecystitis, and cholangitis.